PMDA approval: Halaven Injection 1 mg
Applicant: Eisai Co., Ltd. Approval date: 2016-02-29. Type: Partial change. Active ingredient(s): Eribulin mesylate.
Notes: A drug with a new additional indication for the treatment of soft tissue sarcoma. [Orphan drug]